PMDA approval: Venoglobulin IH 5% I.V. 0.5 g/10 mL Venoglobulin IH 5% I.V. 1 g/20 mL Venoglobulin IH 5% I.V. 2.5 g/50 mL Venoglobulin IH 5% I.V. 5 g/100 mL
Applicant: Benesis Corporation. Approval date: 2011-09-26. Type: Partial change. Active ingredient(s): Polyethylene glycol treated human normal immunoglobulin.
Notes: Drugs with a new additional indication for the treatment of generalized myasthenia gravis (for use only in patients who have not sufficiently responded to steroids or other immunosuppressants). [Orphan drug]